PMDA approval: Padcev for I.V. infusion 20 mg Padcev for I.V. infusion 30 mg
Applicant: Astellas Pharma Inc. Approval date: 2024-09-24. Type: Partial change. Active ingredient(s): Enfortumab vedotin (genetical recombination).
Notes: Drugs with a new indication and a new dosage for the treatment of unresectable urothelial carcinoma. [Priority review]